PMDA approval: Imunace 35 for Injection
Applicant: Shionogi & Co., Ltd. Approval date: 2021-06-23. Type: Partial change. Active ingredient(s): Teceleukin (genetical recombination).
Notes: A drug with a new indication and a new dosage for the enhancement of an antitumor effect of dinutuximab (genetical recombination) for neuroblastoma. [Expedited review]